PMDA approval: Famvir Tab. 250 mg
Applicant: Asahi Kasei Pharma Corporation. Approval date: 2019-02-21. Type: Partial change. Active ingredient(s): Famciclovir.
Notes: A drug with a new dosage indicated for the treatment of herpes simplex.